PMDA approval: Ilaris for S.C. Injection 150 mg
Applicant: Novartis Pharma K.K. Approval date: 2011-09-26. Type: New approval. Active ingredient(s): Canakinumab (genetical recombination).
Notes: A drug with a new active ingredient indicated for the treatment of cryopyrin-associated periodic syndrome. [Orphan drug]